PMDA approval: Synagis for Intramuscular Injection 50 mg Synagis for Intramuscular Injection 100 mg Synagis for Intramuscular Solution 50 mg Synagis for Intramuscular Solution 100 mg
Applicant: Abbvie G.K. Approval date: 2013-08-20. Type: Partial change. Active ingredient(s): Palivizumab (genetical recombination).
Notes: Drugs with new indications for the suppression of development of serious lower respiratory tract disease caused by RS viral infection in newborns, infants, and children aged 24 months or less with immunodeficiency and Down syndrome. [Priority review]